PMDA approval: Comirnaty intramuscular injection
Applicant: Pfizer Japan Inc. Approval date: 2021-02-14. Type: New approval. Active ingredient(s): Tozinameran.
Notes: A drug with a new active ingredient indicated for the prevention of disease caused by SARS-CoV-2 infection (COVID-19). [Drug pertaining to Special Approval for Emergency]